PMDA approval: Herceptin Intravenous Infusion 60 Herceptin Intravenous Infusion 150
Applicant: Chugai Pharmaceutical Co., Ltd. Approval date: 2021-11-25. Type: Partial change. Active ingredient(s): Trastuzumab (genetical recombination).
Notes: Drugs with a new indication and a new dosage for the treatment of unresectable advanced or recurrent HER2-positive salivary gland tumors. [Orphan drug]